PMDA approval: Popscaine 0.5% Inj. 50 mg/10 mL Popscaine 0.5% Inj. syringe 50 mg/10 mL Popscaine 0.25% Inj. 25 mg/10 mL Popscaine 0.25% Inj. syringe 25 mg/10 mL
Applicant: Maruishi Pharmaceutical Co., Ltd. Approval date: 2011-04-22. Type: New approval. Active ingredient(s): Levobupivacaine hydrochloride.
Notes: Drugs with a new route of administration, a new additional indication and new dosages indicated for conduction anesthesia.